PMDA approval: (1) Olumiant Tablets 2 mg (2) Olumiant Tablets 4 mg (3) Olumiant Tablets 1 mg
Applicant: Eli Lilly Japan K.K. Approval date: 2024-03-26. Type: Partial change. Active ingredient(s): Baricitinib (1), (2).
Notes: Drugs with a new additional pediatric dosage and (3) a drug with a new additional pediatric dosage in an additional dosage form, indicated for the treatment of atopic dermatitis in patients who have not responded sufficiently to conventional treatments.